PMDA approval: Zalutia 2.5 mg Tablets Zalutia 5 mg Tablets
Applicant: Eli Lilly Japan K.K. Approval date: 2014-01-17. Type: New approval. Active ingredient(s): Tadalafil.
Notes: Drugs with a new indication and a new dosage in a new additional dosage form indicated for the treatment of dysuria associated with benign prostatic hypertrophy.